PMDA approval: Lynparza Tablets 100 mg Lynparza Tablets 150 mg
Applicant: AstraZeneca K.K. Approval date: 2020-12-25. Type: Partial change. Active ingredient(s): Olaparib.
Notes: Drugs with a new indication and a new dosage for maintenance therapy of unresectable BRCA mutation-positive pancreatic cancer after chemotherapy including treatment with platinum-based antineoplastic drugs. [Orphan drug]